PMDA approval: Unisia Combination Tablets LD Unisia Combination Tablets HD
Applicant: Takeda Pharmaceutical Company Limited. Approval date: 2010-04-16. Type: New approval. Active ingredient(s): Candesartan cilexetil/amlodipine besilate.
Notes: New combination drugs indicated for the treatment of hypertension.